PMDA approval: Fentos Tape 1 mg Fentos Tape 2 mg Fentos Tape 4 mg Fentos Tape 6 mg Fentos Tape 8 mg
Applicant: Hisamitsu Pharmaceutical Co., Inc. Approval date: 2010-04-16. Type: New approval. Active ingredient(s): Fentanyl citrate.
Notes: Drugs with a new route of administration indicated for analgesia in various types of cancer associated with moderate to severe pain which cannot be managed by treatments with non-opioid analgesics or weak opioid analgesics (for use only in patients who switch from an opioid analgesic).